PMDA approval: Cravit Tablets 250 mg Cravit Tablets 500 mg
Applicant: Daiichi Sankyo Co., Ltd. Approval date: 2009-04-22. Type: New approval. Active ingredient(s): Levofloxacin hydrate.
Notes: Drugs in an additional dosage form and with a revised dosage of once-daily administration for conventional indications. 33 Cravit Fine Granules 10% (Daiichi Sankyo Co., Ltd.) Approval